PMDA approval: Botox Vista Injection 50 Units
Applicant: Allergan Japan K.K. Approval date: 2016-05-23. Type: Partial change. Active ingredient(s): Botulinum toxin type.
Notes: A A drug with a new additional indication and a new dosage for the temporary improvement in the appearance of lateral canthal lines (crow's feet lines) in adults aged under 65 years.